PMDA approval: Eylea Intravitreal Injection 40 mg/mL Eylea Intravitreal Injection Kit 40 mg/mL
Applicant: Bayer Yakuhin, Ltd. Approval date: 2015-06-26. Type: Partial change. Active ingredient(s): Aflibercept (genetical recombination).
Notes: Drugs with a revised indication for the treatment of macular edema following retinal vein occlusion.